PMDA approval: Thymoglobuline for Intravenous Infusion 25 mg
Applicant: Sanofi K.K. Approval date: 2014-09-19. Type: Partial change. Active ingredient(s): Anti-human thymocyte immunoglobulin, rabbit.
Notes: A drug with a new additional indication and a new dosage for the treatment of acute rejection after the liver, heart, lungs, pancreas, or small intestinal transplantation.